PMDA approval: Opdivo Intravenous Infusion 20 mg Opdivo Intravenous Infusion 100 mg
Applicant: Ono Pharmaceutical Co., Ltd. Approval date: 2018-05-25. Type: Partial change. Active ingredient(s): Nivolumab (genetical recombination).
Notes: Drugs with a new dosage indicated for the treatment of unresectable melanoma. [Orphan drug]